PMDA approval: Doxil Injection 20 mg
Applicant: Janssen Pharmaceutical K.K. Approval date: 2009-04-22. Type: Partial change. Active ingredient(s): Doxorubicin hydrochloride.
Notes: A drug with a new indication and a new dosage for the treatment of ovarian cancer which has progressed after cancer chemotherapy. [Expedited review]